PMDA approval: Lialda Tab. 1200 mg
Applicant: Mochida Pharmaceutical Co., Ltd. Approval date: 2016-09-28. Type: New approval. Active ingredient(s): Mesalazine.
Notes: A drug in a new dosage form indicated for the treatment of ulcerative colitis (excluding severe cases).